PMDA approval: Beova Tablets 50 mg
Applicant: Kyorin Pharmaceutical Co., Ltd. Approval date: 2018-09-21. Type: New approval. Active ingredient(s): Vibegron.
Notes: A drug with a new active ingredient indicated for the treatment of urinary urgency, urinary frequency, and urge urinary incontinence associated with overactive bladder.